PMDA approval: Cubicin IV 350 mg
Applicant: MSD K.K. Approval date: 2022-06-20. Type: Partial change. Active ingredient(s): Daptomycin.
Notes: A drug with a new additional pediatric dosage indicated for the treatment of sepsis, deep skin infection, secondary infection of trauma, burn, and surgical wounds and secondary infection of erosion and ulcer caused by daptomycin-sensitive methicillin-resistant Staphylococcus aureus (MRSA).